PMDA approval: Ibtrozi Capsules 200 mg
Applicant: Nippon Kayaku Co., Ltd. Approval date: 2025-09-19. Type: New approval. Active ingredient(s): Taletrectinib adipate.
Notes: A drug with a new active ingredient indicated for the treatment of ROS1 fusion gene-positive unresectable advanced or recurrent non-small-cell lung cancer. [Orphan drug]